PMDA approval: Fulmist Intranasal Spray
Applicant: Daiichi Sankyo Company, Limited. Approval date: 2023-03-27. Type: New approval. Active ingredient(s): Live attenuated influenza virus (type A and type B).
Notes: A drug with a new active ingredient indicated for the prevention of influenza.